PMDA approval: Uptravi Tablets 0.2 mg Uptravi Tablets 0.4 mg
Applicant: Nippon Shinyaku Co., Ltd. Approval date: 2021-08-25. Type: Partial change. Active ingredient(s): Selexipag.
Notes: Drugs with a new indication for the treatment of persistent/recurrent chronic thromboembolic pulmonary hypertension (CTEPH) after surgical treatment or inoperable CTEPH. [Orphan drug]